PMDA approval: Tepoxx Capsules 200 mg
Applicant: Japan Biotechno Pharma Co., Ltd. Approval date: 2024-12-27. Type: New approval. Active ingredient(s): Tecovirimat hydrate.
Notes: A drug with a new active ingredient indicated for the treatment of smallpox, mpox, cowpox, as well as complications due to replication of vaccinia virus following vaccination against smallpox. [Expedited review]